PMDA approval: Hydrea Capsules 500 mg
Applicant: Bristol-Myers K.K. Approval date: 2013-03-25. Type: Partial change. Active ingredient(s): Hydroxycarbamide.
Notes: A drug with new additional indications for the treatment of essential thrombocythemia and polycythemia vera. [Public knowledge-based application after PAFSC's preliminary assessment]